PMDA approval: Soliris for Intravenous Infusion 300 mg
Applicant: Alexion Pharma GK. Approval date: 2019-11-22. Type: Partial change. Active ingredient(s): Eculizumab (genetical recombination).
Notes: A drug with a new additional indication for the prevention of relapse of neuromyelitis optica spectrum disorder (including neuromyelitis optica). [Orphan drug]